PMDA approval: Alvesco 50 µg Inhaler 112 puffs Alvesco 100 µg Inhaler 112 puffs Alvesco 200 µg Inhaler 112 puffs Alvesco 200 µg Inhaler 56 puffs
Applicant: Teijin Pharma Ltd. Approval date: 2007-04-18. Type: New approval. Active ingredient(s): Ciclesonide.
Notes: Drugs containing a new active ingredient indicated for the treatment of asthma.